PMDA approval: Venclexta Tablets 10 mg Venclexta Tablets 50 mg Venclexta Tablets 100 mg
Applicant: AbbVie GK. Approval date: 2021-03-23. Type: Partial change. Active ingredient(s): Venetoclax.
Notes: Drugs with a new indication and a new dosage for the treatment of acute myeloid leukemia. [Orphan drug]